PMDA approval: Glivec Tablets 100 mg
Applicant: Novartis Pharma K.K. Approval date: 2012-02-22. Type: Partial change. Active ingredient(s): Imatinib mesilate.
Notes: A drug with new additional indications and a new dosage for the treatment of FIP1L1-PDGFRα- positive hypereosinophilic syndrome or chronic eosinophilic leukemia. [Public knowledge-based application after PAFSC's preliminary assessment] [Orphan drug]